PMDA approval: Vanihep Capsules 150 mg
Applicant: MSD K.K. Approval date: 2014-09-26. Type: New approval. Active ingredient(s): Vaniprevir.
Notes: A drug with a new active ingredient indicated for the improvement of viremia in the following patients with serogroup 1 (genotype I [1a] or II [1b]) chronic hepatitis C: Patients with high levels of blood HCV RNA who are treatment-naive, or Patients who are non-responders or relapsed to therapy including interferon. [Priority review]